PMDA approval: Adcetris for Intravenous Drip Infusion 50 mg
Applicant: Takeda Pharmaceutical Company Limited. Approval date: 2023-11-24. Type: Partial change. Active ingredient(s): Brentuximab vedotin (genetical recombination).
Notes: A drug with a new indication and a new dosage for the treatment of relapsed or refractory CD30-positive cutaneous T-cell lymphoma.